PMDA approval: Elplat I.V. Infusion Solution 50 mg Elplat I.V. Infusion Solution 100 mg Elplat I.V. Infusion Solution 200 mg (Yakult Honsha Co., Ltd.) Oxaliplatin i.v. Infusion 50 mg "Sawai" Oxaliplatin i.v. Infusion 100 mg "Sawai" Oxaliplatin i.v. Infusion 200 mg "Sawai" (Sawai Pharmaceutical Co., Ltd.) Oxaliplatin I.V. Infusion Solution 50 mg "NK" Oxaliplatin I.V. Infusion Solution 100 mg "NK" Oxaliplatin I.V. Infusion Solution 200 mg "NK" (Nippon Kayaku Co., Ltd.) Oxaliplatin I.V. Infusion 50 mg "Nipro" Oxaliplatin I.V. Infusion 100 mg "Nipro" Oxaliplatin I.V. Infusion 200 mg "Nipro" (Nipro Corporation) Oxaliplatin I.V. Drip Infusion 50 mg "DSEP" Oxaliplatin I.V. Drip Infusion 100 mg "DSEP" Oxaliplatin I.V. Drip Infusion 200 mg "DSEP"
Applicant: Daiichi Sankyo Espha Co., Ltd. Approval date: 2018-09-21. Type: Partial change. Active ingredient(s): Oxaliplatin.
Notes: Drugs with a new indication and a new dosage for the treatment of small intestine cancer. [Public knowledge-based application after PAFSC's preliminary assessment]